PMDA approval: Forxiga 5 mg Tablets Forxiga 10 mg Tablets
Applicant: AstraZeneca K.K. Approval date: 2020-11-27. Type: Partial change. Active ingredient(s): Dapagliflozin propylene glycolate hydrate.
Notes: Drugs with a new indication and a new dosage for the treatment of chronic heart failure (for use only in patients receiving standard treatment of chronic heart failure).